PMDA approval: Romiplate for S.C. Injection 250 µg
Applicant: Kyowa Kirin Co., Ltd. Approval date: 2024-11-22. Type: Partial change. Active ingredient(s): Romiplostim (genetical recombination).
Notes: A drug with a new additional pediatric dosage indicated for the treatment of chronic idiopathic thrombocytopenic purpura. [Public knowledge-based application after PAFSC's preliminary assessment]